PMDA approval: Ganirest Subcutaneous 0.25 mg Syringe
Applicant: Nippon Organon K.K. Approval date: 2008-07-16. Type: New approval. Active ingredient(s): Ganirelix acetate.
Notes: A drug containing a new active ingredient indicated for prevention of premature ovulation during controlled ovarian stimulation.